PMDA approval: Fabhalta Capsules 200 mg
Applicant: Novartis Pharma K.K. Approval date: 2025-05-19. Type: Partial change. Active ingredient(s): Iptacopan hydrochloride hydrate.
Notes: A drug with a new indication for the treatment of complement 3 glomerulopathy. [Orphan drug]